PMDA approval: Aricept Tablets 3 mg Aricept Tablets 5 mg Aricept Tablets 10 mg Aricept D Tablets 3 mg Aricept D Tablets 5 mg Aricept D Tablets 10 mg Aricept Fine Granules 0.5% Aricept Oral Jelly 3 mg Aricept Oral Jelly 5 mg Aricept Oral Jelly 10 mg Aricept Dry Syrup 1%
Applicant: Eisai Co., Ltd. Approval date: 2014-09-19. Type: Partial change. Active ingredient(s): Donepezil hydrochloride.
Notes: Drugs with a new additional indication and a new dosage for inhibiting progress of dementia symptoms in patients with dementia with Lewy bodies.